PMDA approval: (1) Lialda Tab. 600 mg (2) Lialda Tab. 1200 mg
Applicant: Mochida Pharmaceutical Co., Ltd. Approval date: 2025-06-24. Type: New approval. Active ingredient(s): Mesalazine.
Notes: (1) A drug with a new dosage in an additional dosage form indicated for the treatment of ulcerative colitis (excluding severe cases). (2) A drug with a new dosage indicated for the treatment of ulcerative colitis (excluding severe cases).